PMDA approval: Amitiza Capsules 24 μg
Applicant: Sucampo Pharma. Ltd. Approval date: 2012-06-29. Type: New approval. Active ingredient(s): Lubiprostone.
Notes: A drug with a new active ingredient indicated for the treatment of chronic constipation (excluding constipation due to organic diseases).